PMDA approval: Humira 40 mg for S.C. Injection Syringe 0.8 mL
Applicant: Abbvie G.K. Approval date: 2013-06-14. Type: Partial change. Active ingredient(s): Adalimumab (genetical recombination).
Notes: A drug with a new additional indication and a new dosage for the treatment of moderate to severe ulcerative colitis (for use only in patients who have not responded sufficiently to conventional treatments).